PMDA approval: Livalo Tablets 1 mg Livalo Tablets 2 mg Livalo OD Tablets 1 mg Livalo OD Tablets 2 mg
Applicant: Kowa Company, Ltd. Approval date: 2015-06-26. Type: Partial change. Active ingredient(s): Pitavastatin calcium.
Notes: Drugs with a new additional pediatric dosage indicated for the treatment of familial hypercholesterolemia.